PMDA approval: NexoBrid Gel 5 g
Applicant: Kaken Pharmaceutical Co., Ltd. Approval date: 2022-12-23. Type: New approval. Active ingredient(s): Purified pineapple stem juice.
Notes: A drug with a new active ingredient indicated for the removal of necrotic tissue of deep dermal burn or deep burn. [Orphan drug]